PMDA approval: Lumakras Tablets 120 mg
Applicant: Amgen K.K. Approval date: 2022-01-20. Type: New approval. Active ingredient(s): Sotorasib.
Notes: A drug with a new active ingredient indicated for the treatment of unresectable advanced or recurrent KRAS G12C mutation-positive non-small cell lung cancer that has progressed after cancer chemotherapy. [Orphan drug]